PMDA approval: Vaxzevria Intramuscular Injection
Applicant: AstraZeneca K.K. Approval date: 2021-05-21. Type: New approval. Active ingredient(s): COVID-19 (SARS-CoV-2) vaccine (recombinant chimpanzee adenovirus vector).
Notes: A drug with a new active ingredient indicated for the prevention of disease caused by SARS-CoV-2 infection (COVID-19). [Drug pertaining to Special Approval for Emergency] Vaccines Nov.11, 2021 143 Comirnaty Intramuscular Injection (Pfizer Japan Inc.) Change Tozinameran A drug with a new dosage indicated for the prevention of disease caused by SARS-CoV-2 infection (COVID-19). [Drug pertaining to Special Approval for Emergency]